PMDA approval: Midafresa Injection 0.1%
Applicant: Alfresa Pharma Corporation. Approval date: 2014-09-26. Type: New approval. Active ingredient(s): Midazolam.
Notes: A drug with a new additional indication and a new dosage in an additional dosage form indicated for the treatment of status epilepticus.